PMDA approval: Lynparza Tablets 100 mg Lynparza Tablets 150 mg
Applicant: AstraZeneca K.K. Approval date: 2022-08-24. Type: Partial change. Active ingredient(s): Olaparib.
Notes: Drugs with a new indication and a new dosage for the postoperative adjuvant treatment of BRCA mutation-positive and HER2-negative breast cancer with a high risk of recurrence. [Orphan drug]